PMDA approval: Maviret Combination Tablets
Applicant: AbbVie GK. Approval date: 2017-09-27. Type: New approval. Active ingredient(s): Glecaprevir hydrate/ Pibrentasvir.
Notes: A new combination drug with a new active ingredient indicated for the improvement of viremia in patients with chronic hepatitis C or compensated cirrhosis type C. [Priority review]